PMDA approval: Jyseleca Tablets 100 mg Jyseleca Tablets 200 mg
Applicant: Gilead Sciences K.K. Approval date: 2020-09-25. Type: New approval. Active ingredient(s): Filgotinib maleate.
Notes: Drugs with a new active ingredient indicated for the treatment of rheumatoid arthritis (including the prevention of structural joint damage) in patients who have not responded sufficiently to conventional treatments.